PMDA approval: Revlimid Capsules 2.5 mg Revlimid Capsules 5 mg
Applicant: Celgene K.K. Approval date: 2017-03-02. Type: Partial change. Active ingredient(s): Lenalidomide hydrate.
Notes: Drugs with a new additional indication and a new dosage for the treatment of relapsed or refractory adult T-cell leukemia/lymphoma. [Orphan drug]